PMDA approval: Plavix Tablets 25 mg Plavix Tablets 75 mg
Applicant: Sanofi-Aventis K.K. Approval date: 2007-10-19. Type: Partial change. Active ingredient(s): Clopidogrel sulfate.
Notes: Drugs with a new indication and dosage for the treatment of acute coronary syndrome (unstable angina and non-ST-elevation myocardial infarction) in patients in whom percutaneous coronary intervention (PCI) is considered. [Priority review]